PMDA approval: Opdivo Intravenous Infusion 20 mg Opdivo Intravenous Infusion 100 mg Opdivo Intravenous Infusion 120 mg Opdivo Intravenous Infusion 240 mg
Applicant: Ono Pharmaceutical Co., Ltd. Approval date: 2021-08-25. Type: Partial change. Active ingredient(s): Nivolumab (genetical recombination).
Notes: Drugs with a new dosage indicated for the treatment of unresectable or metastatic renal cell carcinoma.